PMDA approval: Metgluco Tablets 250 mg Metgluco Tablets 500 mg
Applicant: Sumitomo Dainippon Pharma Co., Ltd. Approval date: 2014-08-29. Type: Partial change. Active ingredient(s): Metformin hydrochloride.
Notes: Drugs with a new dosage indicated for the treatment of type 2 diabetes mellitus (for pediatric use).